PMDA approval: Symproic Tablets 0.2 mg
Applicant: Shionogi & Co., Ltd. Approval date: 2017-03-30. Type: New approval. Active ingredient(s): Naldemedine tosilate.
Notes: A drug with a new active ingredient indicated for the treatment of opioid-induced constipation.